PMDA approval: (1) Nesp Injection 5 µg Plastic Syringe Nesp Injection 10 µg Plastic Syringe Nesp Injection 15 µg Plastic Syringe Nesp Injection 20 µg Plastic Syringe Nesp Injection 30 µg Plastic Syringe Nesp Injection 40 µg Plastic Syringe Nesp Injection 60 µg Plastic Syringe Nesp Injection 120 µg Plastic Syringe Nesp Injection 180 µg Plastic Syringe (2) Darbepoetin Alfa Injection 5 μg Syringe [KKF] Darbepoetin Alfa Injection 10 μg Syringe [KKF] Darbepoetin Alfa Injection 15 μg Syringe [KKF] Darbepoetin Alfa Injection 20 μg Syringe [KKF] Darbepoetin Alfa Injection 30 μg Syringe [KKF] Darbepoetin Alfa Injection 40 μg Syringe [KKF] Darbepoetin Alfa Injection 60 μg Syringe [KKF] Darbepoetin Alfa Injection 120 μg Syringe [KKF] Darbepoetin Alfa Injection 180 μg Syringe [KKF]
Applicant: [1] Kyowa Kirin Co., Ltd., [2] Kyowa Kirin Frontier Co., Ltd. Approval date: 2025-06-24. Type: Partial change. Active ingredient(s): Darbepoetin alfa (genetical recombination).
Notes: Drugs with a new indication and a new dosage for the treatment of anemia resulting from the administration of belzutifan.